PMDA approval: Vectibix for Intravenous Infusion 100 mg Vectibix for Intravenous Infusion 400 mg
Applicant: Takeda Pharmaceutical Company Limited. Approval date: 2025-09-19. Type: Partial change. Active ingredient(s): Panitumumab (genetical recombination).
Notes: Drugs with a new indication and a new dosage for the treatment of unresectable advanced or recurrent KRAS G12C mutation-positive colorectal cancer that has progressed after cancer chemotherapy.